PMDA approval: Riona Tab. 250 mg
Applicant: Japan Tobacco Inc. Approval date: 2021-03-23. Type: Partial change. Active ingredient(s): Ferric citrate hydrate.
Notes: A drug with a new indication and a new dosage for the treatment of iron deficiency anemia.